PMDA approval: Faslodex Intramuscular Injection 250 mg
Applicant: AstraZeneca K.K. Approval date: 2011-09-26. Type: New approval. Active ingredient(s): Fulvestrant.
Notes: A drug with a new active ingredient indicated for the treatment of postmenopausal breast cancer.